PMDA approval: Minirinmelt OD Tablets 120 μg Minirinmelt OD Tablets 240 μg
Applicant: Ferring Pharmaceuticals Co., Ltd. Approval date: 2012-03-30. Type: New approval. Active ingredient(s): Desmopressin acetate hydrate.
Notes: Drugs with a new route of administration indicated for the treatment of nocturnal enuresis associated with decreased urine osmolality or urinary specific gravity.